PMDA approval: Elplat I.V. Infusion Solution 50 mg Elplat I.V. Infusion Solution 100 mg Elplat I.V. Infusion Solution 200 mg
Applicant: Yakult Honsha Co., Ltd. Approval date: 2015-03-20. Type: Partial change. Active ingredient(s): Oxaliplatin.
Notes: Drugs with a new additional indication and a new dosage for the treatment of unresectable advanced or recurrent gastric cancer. [Public knowledge-based application after PAFSC's preliminary assessment]